PMDA approval: Eliquis Tablets 2.5 mg Eliquis Tablets 5 mg
Applicant: Bristol-Myers K.K. Approval date: 2012-12-25. Type: New approval. Active ingredient(s): Apixaban.
Notes: Drugs with a new active ingredient indicated for prevention of ischemic stroke and systemic embolism in patients with nonvalvular atrial fibrillation.